PMDA approval: Endoxan for Injection 100 mg Endoxan for Injection 500 mg
Applicant: Shionogi & Co., Ltd. Approval date: 2024-02-09. Type: Partial change. Active ingredient(s): Cyclophosphamide hydrate.
Notes: Drugs with a new indication and a new dosage for the suppression of graft-versus-host disease in hematopoietic stem cell transplantation. [Public knowledge-based application after PAFSC's preliminary assessment]